PMDA approval: Gabapen Tablets 200 mg Gabapen Tablets 300 mg Gabapen Tablets 400 mg Gabapen Syrop 5%
Applicant: Pfizer Japan Inc. Approval date: 2011-07-01. Type: Partial change. Active ingredient(s): Gabapentine.
Notes: Drugs with a new additional pediatric dosage and an additional dosage form indicated for use as an adjunctive therapy with other antiepileptic drugs to treat partial seizures (including secondary generalized seizures) in patients with epilepsy who have not responded sufficiently to other antiepileptic drugs.